PMDA approval: Nopicor Capsules 2.5 μg
Applicant: Toray Medical Co., Ltd. Approval date: 2014-12-26. Type: New approval. Active ingredient(s): Nalfurafine hydrochloride.
Notes: A drug with a new active ingredient indicated for the improvement of pruritus in patients with chronic liver diseases (for use only when conventional therapies are not sufficiently effective).